PMDA approval: Certican Tablets 0.25 mg Certican Tablets 0.5 mg Certican Tablets 0.75 mg
Applicant: Novartis Pharma K.K. Approval date: 2018-02-23. Type: Partial change. Active ingredient(s): Everolimus.
Notes: Drugs with a new additional indication and a new dosage for the inhibition of rejection in liver transplantation.